PMDA approval: Tagrisso Tablets 40 mg Tagrisso Tablets 80 mg
Applicant: AstraZeneca K.K. Approval date: 2025-05-19. Type: Partial change. Active ingredient(s): Osimertinib mesilate.
Notes: Drugs with a new indication for the maintenance treatment for locally-advanced, unresectable epidermal growth factor receptor (EGFR) mutation-positive non-small cell lung cancer following definitive chemoradiation therapy.